PMDA approval: Izcargo for I.V. Infusion 10 mg
Applicant: JCR Pharmaceuticals Co., Ltd. Approval date: 2021-03-23. Type: New approval. Active ingredient(s): Pabinafusp alfa (genetical recombination).
Notes: A drug with a new active ingredient indicated for the treatment of mucopolysaccharidosis type II. [SAKIGAKE review, orphan drug]